PMDA approval: Fentos Tape 0.5 mg Fentos Tape 1 mg Fentos Tape 2 mg Fentos Tape 4 mg Fentos Tape 6 mg Fentos Tape 8 mg
Applicant: Hisamitsu Pharmaceutical Co., Inc. Approval date: 2020-06-29. Type: Partial change. Active ingredient(s): Fentanyl citrate.
Notes: Drugs with a new indication and a new dosage for the analgesia in various types of cancer with moderate to severe pain that cannot be managed with non-opioid analgesics and weak opioid analgesics.